PMDA approval: Mirapex-LA Tablets 0.375 mg Mirapex-LA Tablets 1.5 mg
Applicant: Nippon Boehringer Ingelheim Co., Ltd. Approval date: 2011-04-22. Type: New approval. Active ingredient(s): Pramipexole hydrochloride hydrate.
Notes: Drugs in new dosage forms and with new dosages indicated for the treatment of Parkinson's disease.